PMDA approval: Kisunla Intravenous Infusion 350 mg
Applicant: Eli Lilly Japan K.K. Approval date: 2025-08-25. Type: Partial change. Active ingredient(s): Donanemab (genetical recombination).
Notes: A drug with a new dosage indicated for slowing the progression of mild cognitive impairment and mild dementia due to Alzheimer’s disease.